PMDA approval: Cell Culture-derived Influenza Emulsion HA Vaccine (prototype) for Intramuscular Injection “Kaketsuken”
Applicant: Kaketsuken [The Chemo-Sero-Therapeutic Research Institute]. Approval date: 2015-03-26. Type: New approval. Active ingredient(s): Cell culture-derived influenza emulsion HA vaccines (prototype).
Notes: A drug with a new active ingredient indicated for the prevention of pandemic influenza. [Orphan drug]